PMDA approval: Ranibizumab BS Intravitreal Injection kit 10 mg/mL "Senju"
Applicant: Senju Pharmaceutical Co., Ltd. Approval date: 2021-09-27. Type: New approval. Active ingredient(s): Ranibizumab (genetical recombination) [ranibizumab biosimilar 1].
Notes: Follow-on biologics indicated for the treatment of age-related macular degeneration associated with subfoveal choroidal neovascularization or choroidal neovascularization in patients with pathologic myopia.